PMDA approval: Ifomide for Injection 1 g
Applicant: Shionogi & Co., Ltd. Approval date: 2012-03-21. Type: Partial change. Active ingredient(s): Ifosfamide.
Notes: A drug with a new additional indication and a new dosage for the treatment of malignant lymphoma. [Public knowledge-based application after PAFSC's preliminary assessment]